PMDA approval: Esanbutol Tablets 125 mg Esanbutol Tablets 250 mg (Sandoz K.K.) Ebutol 125 mg Tablets Ebutol 250 mg Tablets
Applicant: Kaken Pharmaceutical Co., Ltd. Approval date: 2011-05-20. Type: Partial change. Active ingredient(s): Ethambutol hydrochloride.
Notes: Drugs with a new additional indication and a new dosage for the treatment of nontuberculous mycobacteriosis including Mycobacterium avium complex (MAC) infection. [Public knowledge-based application after PAFSC's preliminary assessment]